PMDA approval: Clomid Tablets 50 mg
Applicant: Fuji Pharma Co., Ltd. Approval date: 2022-09-16. Type: Partial change. Active ingredient(s): Clomifene Citrate.
Notes: A drug with a new indication and a new dosage for controlled ovarian stimulation in assisted reproductive technology. [Public knowledge-based application after PAFSC's preliminary assessment]